PMDA approval: MyoMIBG-I123 Injection
Applicant: Fujifilm RI Pharma Co., Ltd. Approval date: 2009-11-06. Type: Partial change. Active ingredient(s): 3-iodobenzylguanidine (123I) injection.
Notes: A drug with a new additional indication and a new dosage for diagnosis of neuroblastoma in tumor scintigraphy.